PMDA approval: Kenketsu Venilon-I for Intravenous Injection 500 mg
Applicant: Kaketsuken [The Chemo-Sero-Therapeutic Research Institute]. Approval date: 2010-01-20. Type: Partial change. Active ingredient(s): Freeze-dried sulfonated human normal immunoglobulin.
Notes: A drug with a new additional indication and a new dosage for the improvement of neurological disorder in patients with Churg-Strauss syndrome or allergic granulomatous angiitis (for use only when steroids are not sufficiently effective). [Orphan drug]